PMDA approval: Avastin 100 mg/4 mL Intravenous Infusion Avastin 400 mg/16 mL Intravenous Infusion
Applicant: Chugai Pharmaceutical Co., Ltd. Approval date: 2013-11-22. Type: Partial change. Active ingredient(s): Bevacizumab (genetical recombination).
Notes: Drugs with a new additional indication for the treatment of ovarian cancer.